PMDA approval: Voxzogo for Subcutaneous Injection 0.4 mg Voxzogo for Subcutaneous Injection 0.56 mg Voxzogo for Subcutaneous Injection 1.2 mg
Applicant: BioMarin Pharmaceutical K.K. Approval date: 2022-06-20. Type: New approval. Active ingredient(s): Vosoritide (genetical recombination).
Notes: Drugs with a new active ingredient indicated for the treatment of achondroplasia prior to epiphyseal closure. [Orphan drug]